PMDA approval: G-Lasta Subcutaneous Injection 3.6 mg
Applicant: Kyowa Hakko Kirin Co., Ltd. Approval date: 2014-09-26. Type: New approval. Active ingredient(s): Pegfilgrastim (genetical recombination).
Notes: A drug with a new active ingredient indicated for decreasing the incidence of cancer chemotherapy-induced febrile neutropenia.